PMDA approval: Sunlenca Subcutaneous Injection 463.5 mg Sunlenca Tablets 300 mg
Applicant: Gilead Sciences, Inc. Approval date: 2023-08-01. Type: New approval. Active ingredient(s): Lenacapavir sodium.
Notes: Drugs with a new active ingredient indicated for the treatment of multidrug-resistant HIV-1 infection. [Orphan drug]